PMDA approval: Nesp Injection 10 µg/1 mL Plastic Syringe Nesp Injection 15 µg/1 mL Plastic Syringe Nesp Injection 20 µg/1 mL Plastic Syringe Nesp Injection 30 µg/1 mL Plastic Syringe Nesp Injection 40 µg/1 mL Plastic Syringe Nesp Injection 60 µg/0.6 mL Plastic Syringe Nesp Injection 120 µg/0.6 mL Plastic Syringe Nesp Injection 180 µg/0.9 mL Plastic Syringe
Applicant: Kyowa Hakko Kirin Co., Ltd. Approval date: 2010-04-16. Type: New approval. Active ingredient(s): Darbepoetin alfa (genetical recombination).
Notes: Drugs with a new route of administration and a new indication for the treatment of renal anemia.